PMDA approval: Celecox Tablets 100 mg Celecox Tablets 200 mg
Applicant: Astellas Pharma Inc. Approval date: 2011-12-22. Type: Partial change. Active ingredient(s): Celecoxib.
Notes: Drugs with a new additional indication and a new dosage for the relief of inflammation and pain after operation, trauma, and tooth extraction.